PMDA approval: Anoro Ellipta 7 doses Anoro Ellipta 30 doses
Applicant: GlaxoSmithKline K.K. Approval date: 2014-07-04. Type: New approval. Active ingredient(s): (1) Umeclidinium bromide/ (2) Vilanterol trifenatate.
Notes: New combination drugs with a new active ingredient indicated for the relief of symptoms secondary to airway obstructive disorder in chronic obstructive pulmonary disease (chronic bronchitis, emphysema) (when a combination treatment of an inhaled long-acting anticholinergic and a long-acting beta-2 agonist is needed).